PMDA approval: Vanflyta Tablets 17.7 mg Vanflyta Tablets 26.5 mg
Applicant: Daiichi Sankyo Company, Limited. Approval date: 2019-06-18. Type: New approval. Active ingredient(s): Quizartinib hydrochloride.
Notes: Drugs with a new active ingredient indicated for the treatment of relapsed or refractory FLT3-ITD mutation-positive acute myeloid leukemia. [Orphan drug]